PMDA approval: Retevmo Capsules 40 mg Retevmo Capsules 80 mg
Applicant: Eli Lilly Japan K.K. Approval date: 2024-06-24. Type: Partial change. Active ingredient(s): Selpercatinib.
Notes: Drugs with a new indication and a new dosage for the treatment of RET fusion gene-positive advanced or recurrent solid tumor. [Orphan drug]